PMDA approval: Requip CR Tablets 2 mg Requip CR Tablets 8 mg
Applicant: GlaxoSmithKline K.K. Approval date: 2012-06-29. Type: New approval. Active ingredient(s): Ropinirole hydrochloride.
Notes: Drugs in new dosage forms and with a new dosage. These drugs are indicated for the treatment of Parkinson's disease.